PMDA approval: Cyramza Injection 100 mg Cyramza Injection 500 mg
Applicant: Eli Lilly Japan K.K. Approval date: 2020-11-27. Type: Partial change. Active ingredient(s): Ramucirumab (genetical recombination).
Notes: Drugs with a new dosage indicated for the treatment of unresectable advanced or recurrent non-small cell lung cancer. Drugs with a new dosage indicated for the treatment of unresectable advanced or recurrent gastric cancer, unresectable advanced or recurrent colon or rectal cancer, unresectable advanced or recurrent non-small cell lung cancer, and unresectable hepatocellular carcinoma whose serum AFP level is greater than 400 ng/mL and conditions have progressed after cancer chemotherapy.